PMDA approval: Xeljanz Tablets 5 mg
Applicant: Pfizer Japan Inc. Approval date: 2018-05-25. Type: Partial change. Active ingredient(s): Tofacitinib citrate.
Notes: A drug with a new additional indication and a new dosage for the remission induction and maintenance therapy of moderate to severe ulcerative colitis (for use only in patients who have not sufficiently responded to conventional treatments).